PMDA approval: Comclo Shampoo 0.05%
Applicant: Maruho Co., Ltd. Approval date: 2017-03-30. Type: New approval. Active ingredient(s): Clobetasol propionate.
Notes: A drug in a new dosage form indicated for the treatment of psoriasis vulgaris of the scalp.